PMDA approval: Koselugo Capsules 10 mg Koselugo Capsules 25 mg
Applicant: Alexion Pharma GK. Approval date: 2022-09-26. Type: New approval. Active ingredient(s): Selumetinib sulfate.
Notes: Drugs with a new active ingredient indicated for the treatment of plexiform neurofibroma in patients with neurofibromatosis type 1. [Orphan drug]